PMDA approval: Lenvima Capsule 4 mg Lenvima Capsule 10 mg
Applicant: Eisai Co., Ltd. Approval date: 2015-03-26. Type: New approval. Active ingredient(s): Lenvatinib mesilate.
Notes: Drugs with a new active ingredient indicated for the treatment of unresectable thyroid cancer. [Orphan drug]